PMDA approval: Novolin R FlexPen Novolin R 100 IU/mL Novolin 30R FlexPen Novolin N FlexPen InnoLet 30R
Applicant: Novo Nordisk Pharma Ltd. Approval date: 2011-03-23. Type: New approval. Active ingredient(s): Insulin human (genetical recombination).
Notes: Drugs with a new active ingredient indicated for the treatment of diabetes mellitus in cases where insulin therapy is indicated. However, this application applies only to the change in manufacturing method of the drug substance, and thus formulation, manufacturing method, indications and dosage and administration of the product are the same as the approved product.